PMDA approval: Allergen Extract for Scratch Test - HDM "TORII" 100,000 JAU/mL
Applicant: Torii Pharmaceutical Co., Ltd. Approval date: 2014-12-26. Type: New approval. Active ingredient(s): Dermatophagoides farinae extract 10,000 AU/mL, Dermatophagoides pteronyssinus extract 10,000 AU/mL.
Notes: A drug with new active ingredients indicated for the identification of allergen in patients with allergic diseases.